PMDA approval: Precedex Intravenous Solution 200 µg "Pfizer" Precedex Intravenous Solution 200 µg/50 mL Syringe "Pfizer"
Applicant: Pfizer Japan Inc. Approval date: 2023-02-24. Type: Partial change. Active ingredient(s): Dexmedetomidine hydrochloride.
Notes: Drugs with a new indication and a new dosage for sedation of non-intubated pediatric patients during non-invasive procedures and examinations. [Drugs for specific use]